PMDA approval: Gonalef 75 Gonalef Pen 300 Gonalef Pen 450 Gonalef Pen 900
Applicant: Merck Serono Co., Ltd. Approval date: 2009-07-07. Type: Partial change. Active ingredient(s): Follitropin alfa (genetical recombination).
Notes: Drugs with a new additional indication and a new dosage for induction of ovulation in patients with anovulation and infrequent ovulation associated with hypothalamic-pituitary dysfunction or polycystic ovarian syndrome.